PMDA approval: Rectabul 2 mg Rectal Foam 14 Doses
Applicant: EA Pharma Co., Ltd. Approval date: 2017-09-27. Type: New approval. Active ingredient(s): Budesonide.
Notes: A drug with a new route of administration indicated for the treatment of ulcerative colitis which is not severe.